PMDA approval: Lamictal Tablets 25 mg Lamictal Tablets 100 mg
Applicant: GlaxoSmithKline K.K. Approval date: 2014-08-29. Type: Partial change. Active ingredient(s): Lamotrigine.
Notes: Drugs with a new additional indication and a new dosage for use in the monotherapy for treatment of partial seizures (including secondary generalized seizure) and tonic-clonic seizures in patients with epilepsy.